PMDA approval: Enhertu for Intravenous Drip Infusion 100 mg
Applicant: Daiichi Sankyo Company, Limited. Approval date: 2020-03-25. Type: New approval. Active ingredient(s): Trastuzumab deruxtecan (genetical recombination).
Notes: A drug with a new active ingredient indicated for the treatment of unresectable or recurrent HER2-positive breast cancer in patients who have previously been treated with chemotherapy (for use only if refractory or intolerant to standard therapies). [Conditional early approval]